PMDA approval: Celecox Tablets 100 mg Celecox Tablets 200 mg
Applicant: Astellas Pharma Inc. Approval date: 2009-06-17. Type: Partial change. Active ingredient(s): Celecoxib.
Notes: Drugs with new additional indications for the treatment of lumbago, scapulohumeral periarthritis, cervico-omo-brachial syndrome, and tendinitis/tenosynovitis.